PMDA approval: Nucala for s.c. Injection 100 mg
Applicant: GlaxoSmithKline K.K. Approval date: 2020-03-25. Type: Partial change. Active ingredient(s): Mepolizumab (genetical recombination).
Notes: A drug with a new additional pediatric dosage indicated for the treatment of bronchial asthma (for use only in patients with intractable bronchial asthma whose asthmatic responses are uncontrollable with conventional therapies).